PMDA approval: Solu-Medrol for Intravenous Use 40 mg Solu-Medrol for Intravenous Use 125 mg Solu-Medrol for Intravenous Use 500 mg Solu-Medrol for Intravenous Use 1000 mg
Applicant: Pfizer Japan Inc. Approval date: 2013-03-25. Type: Partial change. Active ingredient(s): Methylprednisolone sodium succinate.
Notes: Drugs with a new additional indication and a new dosage for the treatment of acute exacerbation of multiple sclerosis. [Public knowledge-based application after PAFSC's preliminary assessment]